PMDA approval: Daunomycin 20 mg for Injection
Applicant: Meiji Seika Pharma Co., Ltd. Approval date: 2021-02-24. Type: Partial change. Active ingredient(s): Daunorubicin hydrochloride.
Notes: A drug with a new dosage indicated for the treatment of acute leukemia (including blast crisis of chronic myeloid leukemia). [Public knowledge-based application]